PMDA approval: Spikevax Intramuscular Injection (previously COVID-19 Vaccine Moderna Intramuscular Injection)
Applicant: Takeda Pharmaceutical Company Limited. Approval date: 2021-12-16. Type: Partial change. Active ingredient(s): Elasomeran (former ingredient name: CX- 024414).
Notes: A drug with a new dosage indicated for the prevention of disease caused by SARS-CoV-2 infection (COVID-19). [Drug pertaining to Special Approval for Emergency]